PMDA approval: Revestive 3.8 mg for S.C. Injection
Applicant: Takeda Pharmaceutical Company Limited. Approval date: 2021-06-23. Type: New approval. Active ingredient(s): Teduglutide (genetical recombination).
Notes: A drug with a new active ingredient indicated for the treatment of short bowel syndrome. [Orphan drug]